PMDA approval: Grazyna Tablets 50 mg
Applicant: MSD K.K. Approval date: 2016-09-28. Type: New approval. Active ingredient(s): Grazoprevir hydrate.
Notes: A drug with a new active ingredient indicated for the improvement of viremia in patients with chronic hepatitis C or compensated cirrhosis type C in serogroup 1 (genotype 1). [Priority review]